PMDA approval: (1) Xylocaine Injection Polyamp 0.5 ％ (AstraZeneca K.K.) (2) Lidocaine Hydrochloride Injection 0.5 ％ [Pfizer]
Applicant: Mylan Seiyaku Ltd. Approval date: 2015-12-21. Type: Partial change. Active ingredient(s): (1) Lidocaine hydrochloride monohydrate (2) Lidocaine hydrochloride.
Notes: Drugs with a new additional indication and a new dosage for the intravenous regional anesthesia in upper extremity surgery. [Public knowledge-based application after PAFSC's preliminary assessment]